PMDA approval: Cabometyx Tablets 20 mg Cabometyx Tablets 60 mg
Applicant: Takeda Pharmaceutical Company Limited. Approval date: 2020-03-25. Type: New approval. Active ingredient(s): Cabozantinib malate.
Notes: Drugs with a new active ingredient indicated for the treatment of unresectable or metastatic renal cell carcinoma.